PMDA approval: Darzquro Combination Subcutaneous Injection
Applicant: Janssen Pharmaceutical K.K. Approval date: 2021-03-23. Type: New approval. Active ingredient(s): Daratumumab (genetical recombination)/ Vorhyaluronidase alfa (genetical recombination).
Notes: A new combination drug with a new active ingredient indicated for the treatment of multiple myeloma.